PMDA approval: Seibule Tablets 25 mg Seibule Tablets 50 mg Seibule Tablets 75 mg
Applicant: Sanwa Kagaku Kenkyusho Co., Ltd. Approval date: 2008-12-22. Type: Partial change. Active ingredient(s): Miglitol.
Notes: Drugs with a new indication for the treatment of postprandial hyperglycemia in Type 2 diabetes mellitus (for use only in patients who have not benefitted sufficiently from taking insulin preparations in conjunction with dietary and exercise regimens) and the treatment of postprandial hyperglycemia in Type 1 diabetes mellitus (for use only in patients who have not benefited sufficiently from dietary and exercise regimens or from taking sulfonylurea or insulin preparations in conjunction with dietary and exercise regimens).